PMDA approval: Velcade Injection 3 mg
Applicant: Janssen Pharmaceutical K.K. Approval date: 2012-12-21. Type: Partial change. Active ingredient(s): Bortezomib.
Notes: A drug with a new route of administration indicated for the treatment of multiple myeloma.